PMDA approval: Lumicef Subcutaneous Injection 210 mg Syringe
Applicant: Kyowa Hakko Kirin Co., Ltd. Approval date: 2016-07-04. Type: New approval. Active ingredient(s): Brodalumab (genetical recombination).
Notes: A drug with a new active ingredient indicated for the treatment of plaque psoriasis, psoriatic arthritis, pustular psoriasis, and erythrodermic psoriasis in patients who have not responded sufficiently to conventional therapies.